PMDA approval: Opdivo Intravenous Infusion 20 mg Opdivo Intravenous Infusion 100 mg Opdivo Intravenous Infusion 240 mg
Applicant: Ono Pharmaceutical Co., Ltd. Approval date: 2020-02-21. Type: Partial change. Active ingredient(s): Nivolumab (genetical recombination).
Notes: 1) Drugs with a new indication for the treatment of unresectable advanced or recurrent microsatellite instability-high (MSI-High) colorectal cancer that has progressed after cancer chemotherapy. 2) Drugs with a new indication for the treatment of unresectable advanced or recurrent oesophageal cancer that has progressed after cancer chemotherapy. 2) [Priority review]